PMDA approval: Relumina Tablets 40 mg
Applicant: Takeda Pharmaceutical Company Limited. Approval date: 2019-01-08. Type: New approval. Active ingredient(s): Relugolix.
Notes: A drug with a new active ingredient indicated for the alleviation of menorrhagia, lower abdominal pain, backache, and anemia that are associated with uterine fibroids.